PMDA approval: Nucala for s.c. Injection 100 mg Syringe Nucala for s.c. Injection 100 mg Pen
Applicant: GlaxoSmithKline K.K. Approval date: 2024-08-28. Type: Partial change. Active ingredient(s): Mepolizumab (genetical recombination).
Notes: Drugs with a new indication and a new dosage for the treatment of chronic rhinosinusitis with nasal polyps (for use only in patients who have not responded sufficiently to conventional treatments).